PMDA approval: Onoact for Intravenous Infusion 50 mg Onoact for Intravenous Infusion 150 mg
Applicant: Ono Pharmaceutical Co., Ltd. Approval date: 2022-08-24. Type: Partial change. Active ingredient(s): Landiolol hydrochloride.
Notes: Drugs with a new indication and a new additional pediatric dosage for the treatment of the following tachyarrhythmia in patients with low cardiac function: supraventricular tachycardia, atrial fibrillation, and atrial flutter.